PMDA approval: Jakavi Tablets 5 mg Jakavi Tablets 10 mg
Applicant: Novartis Pharma K.K. Approval date: 2023-08-23. Type: Partial change. Active ingredient(s): Ruxolitinib phosphate.
Notes: Drugs with a new indication and a new dosage for the treatment of graft-versus-host disease after hematopoietic stem cell transplantation (for use when steroid drugs are not sufficiently effective). [Orphan drug]